PMDA approval: Cymerin 50 mg Injection Cymerin 100 mg Injection
Applicant: Mitsubishi Tanabe Pharma Corporation. Approval date: 2014-12-18. Type: Partial change. Active ingredient(s): Ranimustine.
Notes: Drugs with a new dosage indicated for the treatment of malignant lymphoma. [Expedited review]